PMDA approval: Metgluco Tablets 250 mg
Applicant: Dainippon Sumitomo Pharma Co., Ltd. Approval date: 2010-01-20. Type: New approval. Active ingredient(s): Metformin hydrochloride.
Notes: A drug with a new dosage exceeding the maximum dosage (750 mg/day) of the conventional formulation indicated for the treatment of type 2 diabetes mellitus (for use only in patients who have not responded sufficiently to either [1] diet and exercise therapies alone or [2] sulfonylurea along with diet and exercise therapies).